PMDA approval: Enbrel 25 mg for S.C. Injection
Applicant: Wyeth K.K. Approval date: 2009-07-07. Type: Partial change. Active ingredient(s): Etanercept (genetical recombination).
Notes: A drug with a new indication and a new dosage for the treatment of polyarticular-course juvenile idiopathic arthritis (for use only in patients who have not sufficiently responded to conventional treatments).